PMDA approval: Dayvigo Tablets 2.5 mg Dayvigo Tablets 5 mg Dayvigo Tablets 10 mg
Applicant: Eisai Co., Ltd. Approval date: 2020-01-23. Type: New approval. Active ingredient(s): Lemborexant.
Notes: Drugs with a new active ingredient indicated for the treatment of insomnia.